PMDA approval: Lexapro Tablets 10 mg
Applicant: Mochida Pharmaceutical Co., Ltd. Approval date: 2015-11-20. Type: Partial change. Active ingredient(s): Escitalopram oxalate.
Notes: A drug with a new additional indication for the treatment of social anxiety disorder.